PMDA approval: Jeselhy Tablets 40 mg
Applicant: Taiho Pharmaceutical Co., Ltd. Approval date: 2022-06-20. Type: New approval. Active ingredient(s): Pimitespib.
Notes: A drug with a new active ingredient indicated for the treatment of gastrointestinal stromal tumor that has progressed after cancer chemotherapy.